PMDA approval: Luteum Vaginal Suppository 400 mg
Applicant: Aska Pharmaceutical Co., Ltd. Approval date: 2016-03-28. Type: New approval. Active ingredient(s): Progesterone.
Notes: A drug with a new route of administration indicated for luteal support as part of assisted reproductive technology for infertile women.